PMDA approval: Nexium Capsules 10 mg Nexium Capsules 20 mg Nexium Granules for Suspension 10 mg Nexium Granules for Suspension 20 mg
Applicant: AstraZeneca K.K. Approval date: 2018-01-19. Type: Partial change. Active ingredient(s): Esomeprazole magnesium hydrate.
Notes: Drugs with a new additional pediatric dosage and in additional dosage forms indicated for the treatment of gastric ulcer, duodenal ulcer, anastomotic ulcer, reflux esophagitis, non-erosive reflux disease, and Zollinger-Ellison syndrome.